PMDA approval: Rinvoq Tablets 7.5 mg Rinvoq Tablets 15 mg
Applicant: AbbVie GK. Approval date: 2021-08-25. Type: Partial change. Active ingredient(s): Upadacitinib hydrate.
Notes: Drugs with a new indication and a new dosage for the treatment of atopic dermatitis in patients who have not responded sufficiently to conventional treatments.